PMDA approval: Novastan HI Inj. 10 mg/2 mL (Mitsubishi Tanabe Pharma Corporation) Slonnon HI Injection 10 mg/2 mL
Applicant: Daiichi Sankyo Company, Limited. Approval date: 2011-05-20. Type: Partial change. Active ingredient(s): Argatroban hydrate.
Notes: Drugs with new additional indications and new dosages for the prevention of perfusion blood coagulation during blood extracorporeal circulation (hemodialysis) in patients with heparin-induced thrombocytopenia (HIT) type II and in addition for the prevention of blood coagulation during percutaneous coronary intervention in HIT type II patients (including the patients who have the risk for HIT). [Orphan drug]